PMDA approval: Simulect I.V. Injection 10 mg for Pediatric
Applicant: Novartis Pharma K.K. Approval date: 2008-06-06. Type: Partial change. Active ingredient(s): Basiliximab (genetical recombination).
Notes: A drug with a new dosage and in a new dosage form indicated for supression of acute organ rejection after renal transplantation. [Orphan drug]